PMDA approval: (1) Cravit Tablets 250 mg (2) Cravit Tablets 500 mg (3) Cravit Fine Granules 10％ (Daiichi Sankyo Company, Limited) (4) Levofloxacin Tablets 250 mg “DSEP” (5) Levofloxacin Tablets 500 mg “DSEP” (6) Levofloxacin Fine Granules 10 ％“DSEP”
Applicant: Daiichi Sankyo Espha Co., Ltd. Approval date: 2015-08-24. Type: Partial change. Active ingredient(s): Levofloxacin hydrate.
Notes: Drugs with new additional indications and a new dosage for the treatment of pulmonary tuberculosis and other tuberculosis.